PMDA approval: Alvesco 50 μg Inhaler 112 puffs Alvesco 100 μg Inhaler 112 puffs Alvesco 100 μg Inhaler 56 puffs Alvesco 200 μg Inhaler 56 puffs
Applicant: Teijin Pharma Limited. Approval date: 2011-01-21. Type: Partial change. Active ingredient(s): Ciclesonide.
Notes: Drugs with a new additional pediatric dosage indicated for the treatment of bronchial asthma.